PMDA approval: F-meno Capsules 100 mg
Applicant: Fuji Pharma Co., Ltd. Approval date: 2021-09-27. Type: New approval. Active ingredient(s): Progesterone.
Notes: A drug with a new route of administration indicated for the prevention of endometrial hyperplasia when estrogen preparations were administered for climacteric disturbance or ovarian deficiency symptoms.